PMDA approval: Modiodal Tablets 100 mg
Applicant: Alfresa Pharma Corporation. Approval date: 2011-11-25. Type: Partial change. Active ingredient(s): Modafinil.
Notes: A drug with a new additional indication for the treatment of excessive daytime sleepiness in patients with obstructive sleep apnea syndrome who receive treatment of airway obstruction with continuous positive airway pressure (CPAP) therapy, etc.